PMDA approval: Xeloda Tablets 300
Applicant: Chugai Pharmaceutical Co., Ltd. Approval date: 2020-11-27. Type: Partial change. Active ingredient(s): Capecitabine.
Notes: A drug with a new dosage indicated for the treatment of unresectable or recurrent breast cancer and colorectal cancer. [Public knowledge-based application]